PMDA approval: Calquence Tablets 100 mg
Applicant: AstraZeneca K.K. Approval date: 2024-12-27. Type: New approval. Active ingredient(s): Acalabrutinib maleate hydrate.
Notes: A drug with a new active ingredient indicated for the treatment of chronic lymphocytic leukemia (including small lymphocytic lymphoma).